PMDA approval: Renivace Tablets 2.5 Renivace Tablets 5 Renivace Tablets 10
Applicant: MSD K.K. Approval date: 2012-06-22. Type: Partial change. Active ingredient(s): Enalapril maleate.
Notes: Drugs with a new additional pediatric dosage. These drugs are indicated for the treatment of hypertension. [Public knowledge-based application after PAFSC's preliminary assessment]